PMDA approval: Torisel Injection 25 mg
Applicant: Pfizer Japan Inc. Approval date: 2010-07-23. Type: New approval. Active ingredient(s): Temsirolimus.
Notes: A drug with a new active ingredient indicated for the treatment of unresectable or metastatic renal cell carcinoma.